PMDA approval: Kenketsu Venilon-I for Intravenous Injection 2500 mg
Applicant: Kaketsuken [The Chemo-Sero-Therapeutic Research Institute]. Approval date: 2010-05-13. Type: Partial change. Active ingredient(s): Freeze-dried sulfonated human normal immunoglobulin.
Notes: A drug with a new dosage indicated for the treatment of hypogammaglobulinemia or agammaglobulinemia.